PMDA approval: Adalimumab BS Subcutaneous Injection 40 mg Syringe 0.8 mL “FKB” Adalimumab BS Subcutaneous Injection 40 mg Pen 0.8 mL “FKB” Adalimumab BS Subcutaneous Injection 20 mg Syringe 0.4 mL “FKB”
Applicant: Fujifilm Kyowa Kirin Biologics Co., Ltd. Approval date: 2020-06-29. Type: New approval. Active ingredient(s): Adalimumab (genetical recombination) [adalimumab biosimilar 1].
Notes: Follow-on biologics indicated for the treatment of rheumatoid arthritis (including the prevention of structural joint damage), and the treatment of the following diseases in patients who have not responded sufficiently to conventional treatments; polyarticular-course juvenile idiopathic arthritis, plaque psoriasis, psoriatic arthritis, pustular psoriasis, ankylosing spondylitis and intestinal Behcet’s disease; and the remission induction therapy and maintenance therapy for moderate to severe active Crohn's disease (for use only in patients who have not sufficiently responded to conventional therapies).